PMDA approval: 5-FU Injection 250 mg 5-FU Injection 1000 mg
Applicant: Kyowa Hakko Kirin Co., Ltd. Approval date: 2013-12-20. Type: Partial change. Active ingredient(s): Fluorouracil.
Notes: Drugs with a new additional indication and a new dosage for the treatment of unresectable pancreatic cancer. [Priority review], [Expedited review]